PMDA approval: Cosentyx for S.C. Injection 150 mg Syringe Cosentyx for S.C. Injection 150 mg
Applicant: Novartis Pharma K.K. Approval date: 2014-12-26. Type: New approval. Active ingredient(s): Secukinumab (genetical recombination).
Notes: Drugs with a new active ingredient indicated for the treatment of plaque psoriasis and psoriatic arthritis in patients who have not responded sufficiently to conventional therapies.